PMDA approval: Zosyn for Intravenous Injection 2.25 Zosyn for Intravenous Injection 4.5 Zosyn for I.V. Infusion bag 4.5
Applicant: Taiho Pharmaceutical Co., Ltd. Approval date: 2017-05-18. Type: Partial change. Active ingredient(s): Tazobactam/Piperacillin hydrate.
Notes: Drugs with additional indications and new dosages for the treatment of "bacterial skin infections mainly involving the dermis and/or subcutaneous tissues" and "secondary bacterial infections of pre-existing skin ulcers and/or erosion".